PMDA approval: (1) Fasenra Subcutaneous Injection 30 mg Syringe (2) Fasenra Subcutaneous Injection 10 mg Syringe
Applicant: AstraZeneca K.K. Approval date: 2024-03-26. Type: Partial change. Active ingredient(s): Benralizumab (genetical recombination).
Notes: (1) A drug with a new additional pediatric dosage and (2) a drug with a new additional pediatric dosage in an additional dosage form, indicated for the treatment of bronchial asthma (for use only in patients with intractable bronchial asthma whose asthmatic responses are uncontrollable with conventional therapies).